PMDA approval: Adcirca Tablets 20 mg
Applicant: Eli Lilly Japan K.K. Approval date: 2009-10-16. Type: New approval. Active ingredient(s): Tadalafil.
Notes: A drug with a new indication and a new dosage for the treatment of pulmonary arterial hypertension.